PMDA approval: Darzquro Combination Subcutaneous Injection
Applicant: Janssen Pharmaceutical K.K. Approval date: 2021-08-25. Type: Partial change. Active ingredient(s): Daratumumab (genetical recombination)/ Vorhyaluronidase alfa (genetical recombination).
Notes: A drug with a new indication and a new dosage for the treatment of systemic AL amyloidosis. [Orphan drug]